PMDA approval: TachoSil Tissue Sealing Sheet
Applicant: CSL Behring K.K. Approval date: 2011-09-26. Type: New approval. Active ingredient(s): Human fibrinogen/thrombin fraction.
Notes: A new combination drug indicated for the adhesion and closure of wounds in tissues at surgery in the fields of hepatic surgery, pulmonary surgery, cardiovascular surgery, obstetrics and gynecology, and urologic surgery.